PMDA approval: (1) Estrana Tape 0.72 mg (2) Estrana Tape 0.36 mg
Applicant: Hisamitsu Pharmaceutical Co., Inc. Approval date: 2022-03-11. Type: Partial change. Active ingredient(s): Estradiol.
Notes: (1) A drug with a new indication and a new dosage for adjusting the start timing of controlled ovarian stimulation in assisted reproductive technology and hormone replacement cycle in frozen-thawed embryo transfer. (2) A drug with a new indication and a new dosage for hormone replacement cycle in frozen-thawed embryo transfer. [Public knowledge-based application]